PMDA approval: Besponsa Injection 1 mg
Applicant: Pfizer Japan Inc. Approval date: 2024-03-26. Type: Partial change. Active ingredient(s): Inotuzumab ozogamicin (genetical recombination).
Notes: A drug with a new additional pediatric dosage indicated for the treatment of relapsed or refractory CD22-positive acute lymphoblastic leukemia.